PMDA approval: Fabhalta Capsules 200 mg
Applicant: Novartis Pharma K.K. Approval date: 2024-06-24. Type: New approval. Active ingredient(s): Iptacopan hydrochloride hydrate.
Notes: A drug with a new active ingredient indicated for the treatment of paroxysmal nocturnal haemoglobinuria.